PMDA approval: Gracevit Tablets 50 mg Gracevit Fine Granules 10%
Applicant: Daiichi Sankyo Co., Ltd. Approval date: 2008-01-25. Type: New approval. Active ingredient(s): Sitafloxacin hydrate.
Notes: A drug containing a new active ingredient indicated for the treatment of laryngopharyngitis, tonsillitis (including peritonsillitis and peritonsillar abscess), acute bronchitis, pneumonia, infections secondary to chronic respiratory disease, cystitis, pyelonephritis, urethritis, cervicitis, otitis media, sinusitis, periodontal inflammation, pericoronitis and jaw inflammation.